PMDA approval: Dovato Combination Tablets
Applicant: ViiV Healthcare K.K. Approval date: 2020-01-14. Type: New approval. Active ingredient(s): Dolutegravir sodium/Lamivudine.
Notes: A new combination drug indicated for the treatment of HIV infection. [Orphan drug]